PMDA approval: Mekinist Tablets 0.5 mg Mekinist Tablets 2 mg
Applicant: Novartis Pharma K.K. Approval date: 2018-03-23. Type: Partial change. Active ingredient(s): Trametinib dimethyl sulfoxide.
Notes: Drugs with a new additional indication for the treatment of unresectable advanced or recurrent BRAF mutation-positive non-small-cell lung cancer. [Orphan drug]